PMDA approval: Humira 40 mg for S.C. Injection Syringe 0.8 mL Humira 40 mg for S.C. Injection Syringe 0.4 mL Humira 80 mg for S.C. Injection Syringe 0.8 mL Humira 40 mg for S.C. Injection in pre-filled pen 0.4 mL Humira 80 mg for S.C. Injection in pre-filled pen 0.8 mL
Applicant: AbbVie GK. Approval date: 2018-03-23. Type: Partial change. Active ingredient(s): Adalimumab (genetical recombination).
Notes: Drugs with a new additional indication and a new dosage indicated for the treatment of pustular psoriasis in patients who have not responded sufficiently to conventional therapies.